PMDA approval: Viltepso Intravenous Infusion 250 mg
Applicant: Nippon Shinyaku Co., Ltd. Approval date: 2020-03-25. Type: New approval. Active ingredient(s): Viltolarsen.
Notes: A drug with a new active ingredient indicated for the treatment of Duchenne muscular dystrophy (DMD) with a confirmed deficiency of the dystrophin gene amenable to exon 53 skipping therapy. [SAKIGAKE designation] [Conditional early approval] [Orphan drug]